PMDA approval: Phozevel Tablets 5 mg Phozevel Tablets 10 mg Phozevel Tablets 20 mg Phozevel Tablets 30 mg
Applicant: Kyowa Kirin Co., Ltd. Approval date: 2023-09-25. Type: New approval. Active ingredient(s): Tenapanor hydrochloride.
Notes: Drugs with a new active ingredient indicated for the improvement of hyperphosphatemia in patients with chronic kidney disease on dialysis.